PMDA approval: Enevo Liquid for Enteral Use
Applicant: Abbott Japan Co., Ltd. Approval date: 2014-03-24. Type: New approval. Active ingredient(s): N/A for this combination drug.
Notes: A combination prescription drug with similar formulations indicated for tube feeding for especially patients with long-term oral feeding difficulties. It also generally can be used for nutrient retention for postoperative patients.